PMDA approval: Valtrex Granules 50%
Applicant: GlaxoSmithKline K.K. Approval date: 2007-04-18. Type: Partial change. Active ingredient(s): Valaciclovir hydrochloride.
Notes: A drug with a new indication and dosage for the treatment of varicella in children.